PMDA approval: Avastin 100 mg/4 mL Intravenous Infusion Avastin 400 mg/16 mL Intravenous Infusion
Applicant: Chugai Pharmaceutical Co., Ltd. Approval date: 2020-09-25. Type: Partial change. Active ingredient(s): Bevacizumab (genetical recombination).
Notes: Drugs with a new indication and a new dosage for the treatment of unresectable hepatocellular carcinoma. [Priority review]